PMDA approval: Anafranil Tablets 10 mg Anafranil Tablets 25 mg
Applicant: Alfresa Pharma Corporation. Approval date: 2013-11-22. Type: Partial change. Active ingredient(s): Clomipramine hydrochloride.
Notes: Drugs with a new additional indication and a new dosage for the treatment of cataplexy associated with narcolepsy. [Public knowledge-based application after PAFSC's preliminary assessment]